PMDA approval: Diflucan Capsules 50 mg Diflucan Capsules 100 mg
Applicant: Pfizer Japan Inc. Approval date: 2015-05-26. Type: Partial change. Active ingredient(s): Fluconazole.
Notes: Drugs with new additional indications and a new dosage for the treatment of vaginitis and vulvovaginitis caused by Candida.